PMDA approval: Nesina Tablets 6.25 mg Nesina Tablets 12.5 mg Nesina Tablets 25 mg
Applicant: Takeda Pharmaceutical Company Limited. Approval date: 2010-04-16. Type: New approval. Active ingredient(s): Alogliptin benzoate.
Notes: Drugs with a new active ingredient indicated for the treatment of type 2 diabetes (for use only in patients with inadequate glycemic control by following treatments: 1. Treatment by diet and exercise only 2. In addition to treatment by diet and exercise, treatment by alpha-glucosidase inhibitor).